PMDA approval: Evrysdi Dry Syrup 60 mg
Applicant: Chugai Pharmaceutical Co., Ltd. Approval date: 2021-06-23. Type: New approval. Active ingredient(s): Risdiplam.
Notes: A drug with a new active ingredient indicated for the treatment of spinal muscular atrophy. [Orphan drug]